PMDA approval: Cell Culture-derived Influenza Vaccine (prototype) "Baxter" (Baxter Limited) Cell Culture-derived Influenza Vaccine (prototype) "Takeda" 5 mL
Applicant: Takeda Pharmaceutical Company Limited. Approval date: 2013-04-26. Type: New approval. Active ingredient(s): Cell culture-derived influenza vaccines (prototype).
Notes: Drugs with a new active ingredient indicated for the prevention of pandemic influenza. [Orphan drug]